PMDA approval: Oldamin for Injection 1 g
Applicant: Fuji Chemical Industries Co., Ltd. Approval date: 2017-06-26. Type: Partial change. Active ingredient(s): Monoethanolamine oleate.
Notes: A drug with a new additional indication and a new dosage indicated for the regression of gastric varices.